PMDA approval: Gran Injection 75 Gran Injection 150 Gran Injection M300 Gran Syringe 75 Gran Syringe 150 Gran Syringe M300
Applicant: Kyowa Kirin Co., Ltd. Approval date: 2022-06-20. Type: Partial change. Active ingredient(s): Filgrastim (genetical recombination).
Notes: Drugs with a new indication and a new dosage for use as an adjunctive therapy with antineoplastic agents for the treatment of relapsed or refractory acute myeloid leukemia. [Public knowledge-based application after PAFSC's preliminary assessment]